PMDA approval: Opdivo Intravenous Infusion 20 mg Opdivo Intravenous Infusion 100 mg
Applicant: Ono Pharmaceutical Co., Ltd. Approval date: 2016-12-02. Type: Partial change. Active ingredient(s): Nivolumab (genetical recombination).
Notes: Drugs with a new additional indication for the treatment of relapsed or refractory classical Hodgkin's lymphoma. [Orphan drug]